PMDA approval: (1) Anerem 20 mg for I.V. Injection (2) Anerem 50 mg for I.V. Injection
Applicant: Mundipharma K.K. Approval date: 2025-06-24. Type: New approval. Active ingredient(s): Remimazolam besilate.
Notes: (1) A drug with a new indication and a new dosage in an additional dosage form for sedation during gastrointestinal endoscopy. (2) A drug with a new indication, a new dosage, and other characteristics for sedation during gastrointestinal endoscopy.